PMDA approval: Mektovi Tablets 15 mg
Applicant: Ono Pharmaceutical Co., Ltd. Approval date: 2024-05-17. Type: Partial change. Active ingredient(s): Binimetinib.
Notes: A drug with new indications and a new dosage for the treatments of unresectable BRAF mutation-positive thyroid cancer that has progressed after cancer chemotherapy and unresectable BRAF mutation-positive anaplastic thyroid cancer.